PMDA approval: Lynparza Tablets 100 mg Lynparza Tablets 150 mg
Applicant: AstraZeneca K.K. Approval date: 2019-06-18. Type: Partial change. Active ingredient(s): Olaparib.
Notes: Drugs with a new indication for maintenance treatment of BRCA mutation-positive ovarian cancer in patients who have received first chemotherapy. [Orphan drug]